PMDA approval: Stalevo Combination Tablets L50 Stalevo Combination Tablets L100
Applicant: Novartis Pharma K.K. Approval date: 2014-07-04. Type: New approval. Active ingredient(s): Levodopa/ Carbidopa hydrate/ Entacapone.
Notes: New combination drugs indicated for the treatment of Parkinson's disease [for use in patients having wearing-off phenomenon in the symptoms following administration of levodopa/carbidopa].